PMDA approval: Leustatin Injection 8 mg
Applicant: Janssen Pharmaceutical K.K. Approval date: 2009-03-24. Type: Partial change. Active ingredient(s): Cladribine.
Notes: A drug with a new additional dosage indicated for the treatment of recurrent, relapsing, or refractory indolent B-cell non-Hodgkin's lymphoma including follicular lymphoma, and mantle cell lymphoma.